PMDA approval: Opdivo Intravenous Infusion 20 mg Opdivo Intravenous Infusion 100 mg
Applicant: Ono Pharmaceutical Co., Ltd. Approval date: 2018-08-21. Type: Partial change. Active ingredient(s): Nivolumab (genetical recombination).
Notes: (1) Drugs with new indications and a new dosage for the treatment of unresectable or recurrent malignant pleural mesothelioma and melanoma that have progressed after chemotherapy. [Orphan drug] (2) Drugs with a new dosage indicated for the treatment of melanoma, unresectable or recurrent non-small cell lung cancer, unresectable or metastatic renal cell carcinoma, relapsed or refractory classical Hodgkin lymphoma, recurrent or metastatic head and neck cancer, unresectable or recurrent gastric cancer that has progressed after chemotherapy, and (3) unresectable or metastatic renal cell carcinoma. [(3) Priority review]